PMDA approval: Ranmark Subcutaneous Injection 120 mg
Applicant: Daiichi Sankyo Company, Limited. Approval date: 2014-05-23. Type: Partial change. Active ingredient(s): Denosumab (genetical recombination).
Notes: A drug with a new additional indication and a new dosage for the treatment of giant cell tumor of bone. [Orphan drug]